PMDA approval: Nouriast Tablets 20 mg
Applicant: Kyowa Hakko Kirin Co., Ltd. Approval date: 2013-03-25. Type: New approval. Active ingredient(s): Istradefylline.
Notes: A drug with a new active ingredient indicated for the improvement of wearing-off phenomenon in patients with Parkinson's disease on treatment with levodopa-containing products.